PMDA approval: Ryzodeg Combination Injection FlexTouch Ryzodeg Combination Injection Penfill
Applicant: Novo Nordisk Pharma Ltd. Approval date: 2012-12-25. Type: New approval. Active ingredient(s): Insulin degludec (genetical recombination)/insulin aspart (genetical recombination).
Notes: New combination drugs indicated for the treatment of diabetes mellitus in cases where insulin therapy is indicated.